PMDA approval: Awiqli Injection FlexTouch 300 Units Awiqli Injection FlexTouch 700 Units
Applicant: Novo Nordisk Pharma Ltd. Approval date: 2024-06-24. Type: New approval. Active ingredient(s): Insulin icodec (genetical recombination).
Notes: Drugs with a new active ingredient indicated for the treatment of diabetes mellitus in cases where insulin therapy is indicated.